PMDA approval: Vyndaqel Capsules 20 mg
Applicant: Pfizer Japan Inc. Approval date: 2019-03-26. Type: Partial change. Active ingredient(s): Tafamidis meglumine.
Notes: A drug with a new additional indication and a new dosage for the treatment of transthyretin cardiac amyloidosis (wild-type and hereditary). [SAKIGAKE designation, Orphan drug]